PMDA approval: Litfulo Capsules 50 mg
Applicant: Pfizer Japan Inc. Approval date: 2023-06-26. Type: New approval. Active ingredient(s): Ritlecitinib tosilate.
Notes: A drug with a new active ingredient indicated for the treatment of alopecia areata (for use only in patients with intractable and extensive alopecia areata).